PMDA approval: Abraxane I.V. Infusion 100 mg
Applicant: Taiho Pharmaceutical Co., Ltd. Approval date: 2021-08-25. Type: Partial change. Active ingredient(s): Paclitaxel.
Notes: A drug with a new dosage indicated for the treatment of breast cancer.